PMDA approval: Yervoy Injection 50 mg
Applicant: Bristol-Myers Squibb K.K. Approval date: 2021-05-27. Type: Partial change. Active ingredient(s): Ipilimumab (genetical recombination).
Notes: A drug with a new indication and a new dosage for the treatment of unresectable advanced or recurrent malignant pleural mesothelioma. [Orphan drug]